PMDA approval: Lyrica Capsules 25 mg Lyrica Capsules 75 mg Lyrica Capsules 150 mg
Applicant: Pfizer Japan Inc. Approval date: 2013-02-28. Type: Partial change. Active ingredient(s): Pregabalin.
Notes: Drugs with a new indication for the treatment of neuropathic pain.